PMDA approval: Lazcluze Tablets 80 mg Lazcluze Tablets 240 mg
Applicant: Janssen Pharmaceutical K.K. Approval date: 2025-03-27. Type: New approval. Active ingredient(s): Lazertinib mesilate hydrate.
Notes: Drugs with a new active ingredient indicated for the treatment of unresectable advanced or recurrent epidermal growth factor receptor (EGFR) mutation-positive non-small cell lung cancer.